PMDA approval: Yervoy Injection 50 mg
Applicant: Bristol-Myers Squibb K.K. Approval date: 2018-08-21. Type: Partial change. Active ingredient(s): Ipilimumab (genetical recombination).
Notes: A drug with a new indication and a new dosage for the treatment of unresectable or metastatic renal cell carcinoma. [Priority review]